PMDA approval: Mekinist Tablets 0.5 mg Mekinist Tablets 2 mg
Applicant: Novartis Pharma K.K. Approval date: 2023-11-24. Type: Partial change. Active ingredient(s): Trametinib dimethyl sulfoxide.
Notes: Drugs with a new indication and a new dosage for the treatment of advanced or recurrent BRAF mutation-positive solid tumor (excluding colon/rectal cancer) that is refractory or intolerant to standard therapies and relapsed or refractory BRAF mutation-positive hairy cell leukemia. [Orphan drug]